PMDA approval: Clenafin Topical Solution 10% for Nail
Applicant: Kaken Pharmaceutical Co., Ltd. Approval date: 2014-07-04. Type: New approval. Active ingredient(s): Efinaconazole.
Notes: A drug with a new active ingredient indicated for the treatment of tinea unguium caused by dermatophyte (trichophyton ).